PMDA approval: Kyprolis for Intravenous Injection 10 mg Kyprolis for Intravenous Injection 40 mg
Applicant: Ono Pharmaceutical Co., Ltd. Approval date: 2017-05-18. Type: Partial change. Active ingredient(s): Carfilzomib.
Notes: Drugs with a new dosage and other characteristics indicated for the treatment of relapsed or refractory multiple myeloma. [Orphan drug]